PMDA approval: Apokyn Subcutaneous Injection 30 mg
Applicant: Kyowa Hakko Kirin Co., Ltd. Approval date: 2012-03-30. Type: New approval. Active ingredient(s): Apomorphine hydrochloride hydrate.
Notes: A drug with a new active ingredient indicated for the treatment of "off" episodes associated with Parkinson's disease (when levodopa-containing products that are frequently administered and other anti-Parkinson drugs at increased doses are not sufficiently effective). [Orphan drug]